PMDA approval: Lysakare Injection
Applicant: Fujifilm Toyama Chemical Co., Ltd. Approval date: 2021-06-23. Type: New approval. Active ingredient(s): L-Lysine hydrochloride, L-Arginine hydrochloride.
Notes: A new combination drug indicated for the reduction of renal radiation exposure from Lutetium ( 177Lu) oxodotreotide.